PMDA approval: Imfinzi Injection 120 mg Imfinzi Injection 500 mg
Applicant: AstraZeneca K.K. Approval date: 2018-07-02. Type: New approval. Active ingredient(s): Durvalumab (genetical recombination).
Notes: Drugs with a new active ingredient indicated for the maintenance treatment of locally-advanced, unresectable non-small cell lung cancer following definitive chemoradiation therapy.